PMDA approval: Ultomiris for intravenous Infusion 300 mg
Applicant: Alexion Pharma GK. Approval date: 2020-09-25. Type: Partial change. Active ingredient(s): Ravulizumab (genetical recombination).
Notes: A drug with a new indication and a new dosage for the treatment of atypical hemolytic uremic syndrome (aHUS).